PMDA approval: Sycrest Sublingual Tablets 5 mg Sycrest Sublingual Tablets 10 mg
Applicant: Meiji Seika Pharma Co., Ltd. Approval date: 2016-03-28. Type: New approval. Active ingredient(s): Asenapine maleate.
Notes: Drugs with a new active ingredient indicated for the treatment of schizophrenia.